PMDA approval: Lyrica Capsules 25 mg Lyrica Capsules 75 mg Lyrica Capsules 150 mg
Applicant: Pfizer Japan Inc. Approval date: 2010-10-27. Type: Partial change. Active ingredient(s): Pregabalin.
Notes: Drugs with a new additional indication for the treatment of peripheral neuropathic pain.